PMDA approval: Descovy Combination Tablets LT Descovy Combination Tablets HT
Applicant: Japan Tobacco Inc. Approval date: 2016-12-09. Type: New approval. Active ingredient(s): Emtricitabine/ Tenofovir alafenamide fumarate.
Notes: New combination drugs indicated for the treatment of HIV-1 infection. [Orphan drug]